PMDA approval: Ryjusea Mini ophthalmic solution 0.025%
Applicant: Santen Pharmaceutical Co., Ltd. Approval date: 2024-12-27. Type: New approval. Active ingredient(s): Atropine sulfate hydrate.
Notes: A drug with a new indication in a new dosage form for slowing the progression of myopia.